PMDA approval: Lyumjev Cart Lyumjev MirioPen Lyumjev MirioPen HD Lyumjev Injection 100 units/mL
Applicant: Eli Lilly Japan K.K. Approval date: 2020-03-25. Type: New approval. Active ingredient(s): Insulin lispro (genetical recombination).
Notes: Drugs in new dosage forms indicated for the treatment of diabetes mellitus in cases where insulin therapy is indicated.